PMDA approval: Tabrecta Tablets 150 mg Tabrecta Tablets 200 mg
Applicant: Novartis Pharma K.K. Approval date: 2020-06-29. Type: New approval. Active ingredient(s): Capmatinib hydrochloride hydrate.
Notes: Drugs with a new active ingredient indicated for the treatment of unresectable advanced or recurrent MET exon 14 (METex14) skipping mutation-positive non-small-cell lung cancer. [Orphan drug]